PMDA approval: Xeomin 50 Units for Intramuscular Injection Xeomin 100 Units for Intramuscular Injection Xeomin 200 Units for Intramuscular Injection
Applicant: Teijin Pharma Limited. Approval date: 2021-06-23. Type: Partial change. Active ingredient(s): Incobotulinumtoxin A.
Notes: Drugs with a new indication and a new dosage for the treatment of lower limb spasticity.